PMDA approval: Laserphyrin 100 mg for Injection
Applicant: Meiji Seika Pharma Co., Ltd. Approval date: 2015-05-26. Type: Partial change. Active ingredient(s): Talaporfin sodium.
Notes: A drug with a new additional indication for the treatment of recurrent esophageal cancer associated with local persistence after chemoradiotherapy or radiotherapy. [Orphan drug]